PMDA approval: Tremfya Subcutaneous Injection 100 mg Syringe
Applicant: Janssen Pharmaceutical K.K. Approval date: 2018-03-23. Type: New approval. Active ingredient(s): Guselkumab (genetical recombination).
Notes: A drug with a new active ingredient indicated for the treatment of plaque psoriasis, psoriatic arthritis, pustular psoriasis, and erythrodermic psoriasis in patients who have not responded sufficiently to conventional therapies.